PMDA approval: (1) Rinvoq Tablets 7.5 mg (2) Rinvoq Tablets 15 mg (3) Rinvoq Tablets 30 mg (4) Rinvoq Tablets 45 mg
Applicant: AbbVie GK. Approval date: 2023-06-26. Type: Partial change. Active ingredient(s): Upadacitinib hydrate (1) (2) (3).
Notes: Drugs with a new indication and a new dosage for the remission induction therapy and the maintenance therapy of moderate to severe active Crohn’s disease (for use only in patients who have not sufficiently responded to conventional treatments). (4) A drug with a new indication and a new dosage for the remission induction therapy of moderate to severe active Crohn’s disease (for use only in patients who have not sufficiently responded to conventional treatments).